PMDA approval: Aflibercept BS Solution for Intravitreal Injection 40 mg/mL "SCD" Aflibercept BS Kit for Intravitreal Injection 40 mg/mL "SCD"
Applicant: SamChunDang Pharm. Co., Ltd. Approval date: 2025-09-19. Type: New approval. Active ingredient(s): Aflibercept (genetical recombination) [aflibercept biosimilar 3].
Notes: Biosimilars indicated for the treatment of age-related macular degeneration associated with subfoveal choroidal neovascularization, macular edema following retinal vein occlusion, and choroidal neovascularization in patients with pathologic myopia.